PMDA approval: Axumin Injection
Applicant: Nihon Medi-Physics Co., Ltd. Approval date: 2021-03-23. Type: New approval. Active ingredient(s): Fluciclovine (18F).
Notes: A drug with a new active ingredient indicated for the visualization of tumors in patients with suspected to be newly identified malignant glioma, (for use only in supporting determination of the extent of tumor resection during tumor resection planning using magnetic resonance imaging).